PMDA approval: Meropen for Intravenous Drip Infusion Vial 0.25 g Meropen for Intravenous Drip Infusion Vial 0.5 g Meropen for Intravenous Drip Infusion Kit 0.5 g
Applicant: Dainippon Sumitomo Pharma Co., Ltd. Approval date: 2011-03-10. Type: Partial change. Active ingredient(s): Meropenem hydrate.
Notes: Drugs with a new dosage (the maximum daily dose has been changed) in patients with general infections that are severe and refractory.